PMDA approval: Glash Vista Solution for external use 0.03% 3 mL Glash Vista Solution for external use 0.03% 5 mL
Applicant: Allergan Japan K.K. Approval date: 2014-03-24. Type: New approval. Active ingredient(s): Bimatoprost.
Notes: Drugs with a new route of administration for the treatment of eyelashe hypotrichosis.